PMDA approval: Ilaris Solution for S.C. Injection 150 mg
Applicant: Novartis Pharma K.K. Approval date: 2025-03-27. Type: Partial change. Active ingredient(s): Canakinumab (genetical recombination).
Notes: A drug with a new indication and a new dosage for the treatment of adult-onset Still's disease in patients who have not responded sufficiently to conventional treatments. [Orphan drug]